PMDA approval: Zyprexa for Intramuscular Injection 10 mg
Applicant: Eli Lilly Japan K.K. Approval date: 2012-09-28. Type: New approval. Active ingredient(s): Olanzapine.
Notes: A drug with a new route of administration indicated for the treatment of acute agitation associated with schizophrenia.